PMDA approval: Onecrinone 90mg Progesterone vaginal gel
Applicant: Merck Serono Co., Ltd. Approval date: 2016-07-04. Type: New approval. Active ingredient(s): Progesterone.
Notes: A drug with a new route of administration indicated for luteal support as part of assisted reproductive technology for infertile women.